PMDA approval: Fastic Tablets 30 Fastic Tablets 90 (Ajinomoto Co., Inc.) Starsis Tablets 30 mg Starsis Tablets 90 mg
Applicant: Astellas Pharma Inc. Approval date: 2008-12-22. Type: Partial change. Active ingredient(s): Nateglinide.
Notes: Drugs with a new additional indication for the treatment of postprandial glucose excursions in Type 2 diabetes mellitus (for use only when treatment with thiazolidines in conjunction with dietary and exercise regimens is not sufficiently effective).